PMDA approval: Zolinza Capsules 100 mg
Applicant: MSD K.K. Approval date: 2011-07-01. Type: New approval. Active ingredient(s): Vorinostat.
Notes: A drug with a new active ingredient indicated for the treatment of cutaneous T-cell lymphoma.